PMDA approval: Spiriva 2.5 μg Respimat 60 puffs
Applicant: Nippon Boehringer Ingelheim Co., Ltd. Approval date: 2010-01-20. Type: New approval. Active ingredient(s): Tiotropium bromide hydrate.
Notes: A drug in a new dosage form and with a new dosage as a kit product consisting of Respimat inhaler and a cartridge (solution).